PMDA approval: Actemra 80 mg for Intravenous Infusion Actemra 200 mg for Intravenous Infusion Actemra 400 mg for Intravenous Infusion
Applicant: Chugai Pharmaceutical Co., Ltd. Approval date: 2008-04-16. Type: New approval. Active ingredient(s): Tocilizumab (genetical recombination).
Notes: Drugs with a new indication and a new dosage in a new dosage form for the treatment of rheumatoid arthritis (including prevention of structural damage of joint) that cannot be treated sufficiently with conventional therapies, active juvenile idiopathic arthritis in multiple joints, and systemic juvenile idiopathic arthritis. [Priority review] [Expedited review]